PMDA approval: Insulin Glargine BS Inj. Cartridges [Lilly] Insulin Glargine BS Inj. MirioPen [Lilly]
Applicant: Eli Lilly Japan K.K. Approval date: 2014-12-26. Type: New approval. Active ingredient(s): Insulin glargine (genetical recombination) [Insulin glargine biosimilar 1].
Notes: Follow-on biologics indicated for the treatment of diabetes mellitus where insulin therapy is indicated.